PMDA approval: Samsca Tablets 7.5 mg
Applicant: Otsuka Pharmaceutical Co., Ltd. Approval date: 2013-09-13. Type: Partial change. Active ingredient(s): Tolvaptan.
Notes: A drug with a new additional indication and a new dosage for the treatment of fluid retention in patients with hepatic cirrhosis who have not responded sufficiently to other diuretics such as loop diuretics.